PMDA approval: Allydone Patches 27.5 mg Allydone Patches 55 mg
Applicant: Teikoku Seiyaku Co., Ltd. Approval date: 2022-12-23. Type: New approval. Active ingredient(s): Donepezil.
Notes: Drugs with a new active ingredient indicated for inhibiting progression of symptoms of dementia in patients with Alzheimer's disease.